PMDA approval: Xultophy Combination Injection FlexTouch
Applicant: Novo Nordisk Pharma Ltd. Approval date: 2019-06-18. Type: New approval. Active ingredient(s): Insulin degludec (genetical recombination) /Liraglutide (genetical recombination).
Notes: A new combination drug indicated for the treatment of type 2 diabetes mellitus in cases where insulin therapy is indicated.